PMDA approval: Stelara Subcutaneous Injection 45 mg
Applicant: Janssen Pharmaceutical K.K. Approval date: 2011-01-21. Type: New approval. Active ingredient(s): Ustekinumab (genetical recombination).
Notes: A drug with a new active ingredient indicated for the treatment of plaque psoriasis and psoriatic arthritis in patients who have not responded sufficiently to conventional treatments.